PMDA approval: Claforan Injection 0.5 g Claforan Injection 1 g (Sanofi-Aventis K.K.) Cefotax Injection 0.5 g Cefotax Injection 1 g
Applicant: Sanofi-Aventis Nichi-Iko K.K. Approval date: 2011-12-22. Type: Partial change. Active ingredient(s): Cefotaxime sodium.
Notes: Drugs with a new dosage for the treatment of pediatric purulent meningitis. [Public knowledge-based application after PAFSC's preliminary assessment]